PMDA approval: Minirinmelt OD Tablets 120 μg Minirinmelt OD Tablets 240 μg
Applicant: Ferring Pharmaceuticals Co., Ltd. Approval date: 2012-12-21. Type: Partial change. Active ingredient(s): Desmopressin acetate hydrate.
Notes: Drugs with a new indication and a new dosage for the treatment of central diabetes insipidus.